PMDA approval: Byclot Combination Intravenous Injection
Applicant: KM Biologics Co., Ltd. Approval date: 2022-08-24. Type: Partial change. Active ingredient(s): Freeze-dried activated human blood coagulation factor VII concentrate containing factor X.
Notes: A drug with a new indication and a new dosage for the control of bleeding tendency in patients who have inhibitors against blood coagulation factor VIII or IX.